PMDA approval: Freeze-dried Live Attenuated Varicella Vaccine "Biken"
Applicant: The Research Foundation for Microbial Diseases of Osaka University. Approval date: 2016-03-18. Type: Partial change. Active ingredient(s): Freeze-dried live attenuated varicella vaccine.
Notes: A drug with a new additional indication for the prevention of herpes zoster in individuals 50 years of age and older.